PMDA approval: (1) Vimpat Dry Syrup 10％ (2) Vimpat Tablets 50 mg (3) Vimpat Tablets 100 mg (4) Vimpat for I.V. Infusion 200 mg
Applicant: UCB Japan Co., Ltd. Approval date: 2019-01-08. Type: New approval. Active ingredient(s): Lacosamide (1)-(3).
Notes: A drug in an additional dosage form and drugs with a new additional pediatric dosage indicated for the treatment of partial seizures (including secondary generalized seizures) in patients with epilepsy. (4) A drug with a new route of administration indicated for the treatment of partial seizures (including secondary generalized seizures) in patients with epilepsy. It is used as an alternative therapy for lacosamide oral formulation in patients who are temporarily unable to be administered orally.